PMDA approval: Minclea Catapasm for Internal Use 0.8%
Applicant: Nihon Pharmaceutical Co., Ltd. Approval date: 2010-10-27. Type: New approval. Active ingredient(s): l-Menthol.
Notes: A drug with a new additional indication and a new dosage and in an additional dosage form for the inhibition of gastric peristalsis in endoscopy of the upper gastrointestinal tract.